PMDA approval: Sumiferon 300 Sumiferon 600 Sumiferon DS 300 Sumiferon DS 600
Applicant: Dainippon Sumitomo Pharma Co., Ltd. Approval date: 2008-10-16. Type: Partial change. Active ingredient(s): Interferon alfa (NAMALWA).
Notes: Drugs with a new indication and a new dosage for the treatment of viremia in patients with compensated cirrhosis type C (excluding patients with high blood levels of serogroup 1 HCV-RNA).